PMDA approval: Lucentis Solution for Intravitreal Injection 2.3 mg/0.23 mL
Applicant: Novartis Pharma K.K. Approval date: 2013-08-20. Type: Partial change. Active ingredient(s): Ranibizumab (genetical recombination).
Notes: A drug with new additional indications and a new dosage for the treatment of macular edema following retinal vein occlusion and choroidal neovascularisasion in pathologic myopia.